PMDA approval: Artist Tablets 2.5 mg Artist Tablets 10 mg Artist Tablets 20 mg
Applicant: Daiichi Sankyo Company, Limited. Approval date: 2015-08-24. Type: Partial change. Active ingredient(s): Carvedilol.
Notes: Drugs with a new additional indication and a new dosage for the treatment of tachycardiac atrial fibrillation.